PMDA approval: Spiolto Respimat 28 puffs Spiolto Respimat 60 puffs
Applicant: Nippon Boehringer Ingelheim Co., Ltd. Approval date: 2015-09-28. Type: New approval. Active ingredient(s): Tiotropium bromide hydrate/Olodaterol hydrochloride.
Notes: New combination drugs with a new active ingredient indicated for the relief of symptoms secondary to airway obstructive disorder in chronic obstructive pulmonary disease (chronic bronchitis, emphysema) (when a combination treatment of an inhaled long-acting anticholinergic and a long-acting beta-2 agonist is needed).